PMDA approval: 5-FU Injection 250 mg 5-FU Injection 1000 mg
Applicant: Kyowa Kirin Co., Ltd. Approval date: 2023-03-27. Type: Partial change. Active ingredient(s): Fluorouracil.
Notes: Drugs with a new indication and a new dosage for the treatment of unresectable advanced or recurrent gastric cancer. [Public knowledge-based application after PAFSC's preliminary assessment]